PMDA approval: Teribone Inj. 56.5 μg
Applicant: Asahi Kasei Pharma Corporation. Approval date: 2017-05-18. Type: Partial change. Active ingredient(s): Teriparatide acetate.
Notes: A drug with a new dosage indicated for the treatment of osteoporosis with high risk of fracture.